PMDA approval: Filgrastim BS Inj. 75 μg Syringe "NK" Filgrastim BS Inj. 150 μg Syringe "NK" Filgrastim BS Inj. 300 μg Syringe "NK" (Nippon Kayaku Co., Ltd.) Filgrastim BS Injection 75 μg Syringe "Teva" Filgrastim BS Injection 150 μg Syringe "Teva" Filgrastim BS Injection 300 μg Syringe "Teva"
Applicant: Teva Pharma Japan Inc. Approval date: 2013-02-28. Type: New approval. Active ingredient(s): Filgrastim (genetical recombination) [filgrastim biosimilar 2].
Notes: Follow-on biologics indicated for mobilization of hematopoietic stem cells to peripheral blood, promotion of increases in neutrophil count at the time of hematopoietic stem cell transplantation, and the treatment of neutropenia caused by cancer chemotherapy, neutropenia which affects the treatment of human immunodeficiency virus (HIV) infection, neutropenia associated with myelodysplastic syndrome, neutropenia associated with aplastic anemia, and congenital/idiopathic neutropenia.